PMDA approval: Imfinzi Injection 120 mg Imfinzi Injection 500 mg
Applicant: AstraZeneca K.K. Approval date: 2022-12-23. Type: Partial change. Active ingredient(s): Durvalumab (genetical recombination).
Notes: Drugs with a new indication and a new dosage for the treatment of unresectable biliary tract cancer. [Orphan drug]